PMDA approval: Yaz Combination Tablets
Applicant: Bayer Yakuhin, Ltd. Approval date: 2010-07-23. Type: New approval. Active ingredient(s): Drospirenone/ethinylestradiol betadex.
Notes: A new combination drug with a new active ingredient indicated for the treatment of dysmenorrhea.